PMDA approval: Azimycin Ophthalmic Solution 1%
Applicant: Senju Pharmaceutical Co., Ltd. Approval date: 2019-06-18. Type: New approval. Active ingredient(s): Azithromycin hydrate.
Notes: A drug with a new route of administration indicated for the treatment of conjunctivitis, blepharitis, hordeolum and dacryocystitis caused by azithromycin-sensitive Staphylococcus spp., Streptococcus spp., Streptococcus pneumoniae, Corynebacterium spp., Haemophilus influenzae or Propionibacterium acnes.